PMDA approval: Endoxan for Injection 100 mg Endoxan for Injection 500 mg
Applicant: Shionogi & Co., Ltd. Approval date: 2015-06-26. Type: Partial change. Active ingredient(s): Cyclophosphamide hydrate.
Notes: Drugs with a revised indication and a new dosage for the relief of symptoms of malignant lymphoma. [Expedited review]